PMDA approval: Vepesid Capsules 25 mg Vepesid Capsules 50 mg (Bristol-Myers K.K.) Lastet S Cap. 25 mg Lastet S Cap. 50 mg
Applicant: Nippon Kayaku Co., Ltd. Approval date: 2012-02-22. Type: Partial change. Active ingredient(s): Etoposide.
Notes: Drugs with a new additional indication and a new dosage for the treatment of ovarian cancer which has progressed after cancer chemotherapy. [Public knowledge-based application after PAFSC's preliminary assessment]